PMDA approval: Taurine powder 98% “Taisho”
Applicant: Taisho Pharmaceutical Co., Ltd. Approval date: 2019-02-21. Type: Partial change. Active ingredient(s): Taurine.
Notes: A drug with a new additional indication and a new dosage for the inhibition of stroke-like episodes in patients with mitochondrial myopathy, encephalopathy, lactic acidosis and stroke-like episodes (MELAS). [Orphan drug]